PMDA approval: Influenza HA Vaccine "Biken" Flubik HA Flubik HA Syringe
Applicant: The Research Foundation for Microbial Diseases of Osaka University. Approval date: 2011-08-08. Type: Partial change. Active ingredient(s): Influenza HA Vaccine.
Notes: Drugs with a new pediatric dosage indicated for the prevention of influenza. [Expedited review]